PMDA approval: Pralia Subcutaneous Injection 60 mg Syringe
Applicant: Daiichi Sankyo Company, Limited. Approval date: 2013-03-25. Type: New approval. Active ingredient(s): Denosumab (genetical recombination).
Notes: A drug with a new indication and a new dosage for the treatment of osteoporosis.